PMDA approval: Lanthanum Carbonate Granules 250 mg "Nipro" Lanthanum Carbonate Granules 500 mg "Nipro"
Applicant: Nipro Corporation. Approval date: 2021-03-23. Type: Partial change. Active ingredient(s): Lanthanum carbonate hydrate.
Notes: Drugs with a new indication for the improvement of hyperphosphatemia associated with administration of FGFR inhibitor. [Expedited review]